PMDA approval: Viekirax Combination Tablets
Applicant: AbbVie GK. Approval date: 2016-09-28. Type: Partial change. Active ingredient(s): Ombitasvir hydrate/ Paritaprevir hydrate/ Ritonavir.
Notes: A drug with a new additional indication and a new dosage for the improvement of viremia in patients with chronic hepatitis C in serogroup 2 (genotype 2).